PMDA approval: Elrexfio S.C. Injection 44 mg Elrexfio S.C. Injection 76 mg
Applicant: Pfizer Japan Inc. Approval date: 2025-06-24. Type: Partial change. Active ingredient(s): Elranatamab (genetical recombination).
Notes: Drugs with a new dosage indicated for the treatment of relapsed or refractory multiple myeloma (for use only if refractory or intolerant to standard therapies). [Orphan drug]